PMDA approval: Herceptin Intravenous Infusion 60 Herceptin Intravenous Infusion 150
Applicant: Chugai Pharmaceutical Co., Ltd. Approval date: 2013-06-14. Type: Partial change. Active ingredient(s): Trastuzumab (genetical recombination).
Notes: Drugs with a new dosage for the treatment of breast cancer with HER2 overexpression. [Public knowledge-based application after PAFSC's preliminary assessment]